PMDA approval: Azorga Combination Ophthalmic Suspension
Applicant: Alcon Japan Ltd. Approval date: 2013-09-20. Type: New approval. Active ingredient(s): (1) Brinzolamide (2) Timolol maleate.
Notes: A new combination drug indicated for the treatment of glaucoma and ocular hypertension in patients who have not responded sufficiently to other glaucoma drugs.